PMDA approval: Orphacol 50 mg Capsules
Applicant: ReqMed Company, Ltd. Approval date: 2023-03-27. Type: New approval. Active ingredient(s): Cholic acid.
Notes: A drug with a new active ingredient indicated for the treatment of inborn errors of bile acid metabolism. [Orphan drug]